PMDA approval: Elneopa No. 1 Injection Elneopa No. 2 Injection
Applicant: Otsuka Pharmaceutical Factory, Inc. Approval date: 2009-03-31. Type: New approval. Active ingredient(s): N/A for this combination drug.
Notes: A combination drug similar to other products indicated for the supplementation of water, electrolytes, calories (a calorie source), amino acids, vitamins, Zn, Fe, Mn, and I in patients in whom peroral or enteral nutrition is impossible or insufficient, and who have to rely on parenteral nutrition.